PMDA approval: Sawacillin Fine Granules 10% Sawacillin Capsules 125 Sawacillin Capsules 250 Sawacillin Tablets 250 (Astellas Pharma Inc.) Pasetocin Fine Granules 10% Pasetocin Capsules 125 Pasetocin Capsules 250 Pasetocin Tablets 250
Applicant: Kyowa Hakko Kirin Co., Ltd. Approval date: 2012-02-22. Type: Partial change. Active ingredient(s): Amoxicillin hydrate.
Notes: Drugs with a revised maximum dosage for pediatric patients indicated for the treatment of infections except Helicobacter pylori infection. [Public knowledge-based application after PAFSC's preliminary assessment]